PMDA approval: Levitra Tablets 5 mg Levitra Tablets 10 mg Levitra Tablets 20 mg
Applicant: Bayer Yakuhin Ltd. Approval date: 2007-05-24. Type: Partial change. Active ingredient(s): Vardenafil hydrochloride hydrate.
Notes: Drugs with a new dosage indicated for the treatment of erectile dysfunction (patients unable to obtain or maintain erections sufficient for satisfactory sexual activity).